PMDA approval: Canalia Combination Tablets
Applicant: Mitsubishi Tanabe Pharma Corporation. Approval date: 2017-07-03. Type: New approval. Active ingredient(s): Teneligliptin hydrobromide hydrate/Canagliflozin hydrate.
Notes: A new combination drug indicated for the treatment of type 2 diabetes mellitus (only when a concomitant use of teneligliptin hydrobromide hydrate with canagliflozin hydrate is deemed appropriate).